PMDA approval: Neoral 10 mg Capsules Neoral Solution 10% Neoral 25 mg Capsules Neoral 50 mg Capsules
Applicant: Novartis Pharma K.K. Approval date: 2013-03-25. Type: Partial change. Active ingredient(s): Ciclosporin.
Notes: Drugs with a new indication and a new dosage for the treatment of non-Behcet's, non-infectious uveitis (active non-infectious uveitis in the intermediate or posterior area in patients who have not responded sufficiently to conventional treatments and may involve deterioration of visual acuity). [Public knowledge-based application after PAFSC's preliminary assessment]